PMDA approval: Trelegy 100 Ellipta 14 doses Trelegy 100 Ellipta 30 doses
Applicant: GlaxoSmithKline K.K. Approval date: 2019-03-26. Type: New approval. Active ingredient(s): Fluticasone furoate/Umeclidinium bromide/Vilanterol trifenatate.
Notes: New combination drugs indicated for the relief of symptoms in patients with chronic obstructive pulmonary disease (chronic bronchitis, emphysema) (who require a combination therapy with an inhaled corticosteroid, a long-acting inhaled anticholinergic agent and a long-acting beta-2 agonist).